PMDA approval: Fosrenol Chewable Tablets 250 mg Fosrenol Chewable Tablets 500 mg
Applicant: Bayer Yakuhin Ltd. Approval date: 2008-10-16. Type: New approval. Active ingredient(s): lanthanum carbonate hydrate.
Notes: Drugs containing a new active ingredient indicated for the treatment of hyperphosphatemia in dialysis patients with chronic renal failure.